PMDA approval: Fasenra Subcutaneous Injection 30 mg Syringe
Applicant: AstraZeneca K.K. Approval date: 2018-01-19. Type: New approval. Active ingredient(s): Benralizumab (genetical recombination).
Notes: A drug with a new active ingredient indicated for the treatment of bronchial asthma (for use only in patients with intractable bronchial asthma whose asthmatic responses are uncontrollable with conventional therapies).